PMDA approval: Adsorbed Cell Culture-derived Influenza Vaccine H5N1 for Intramuscular Injection 30μg/mL “Kitasato Daiichi Sankyo”
Applicant: Kitasato Daiichi Sankyo Vaccine Co., Ltd. Approval date: 2016-03-18. Type: Partial change. Active ingredient(s): Adsorbed cell culture-derived influenza vaccine (H5N1).
Notes: A drug with a new additional pediatric dosage indicated for the prevention of pandemic influenza (H5N1). [Orphan drug]